PMDA approval: Nasonex Nasal 50 μg 56 sprays Nasonex Nasal 50 μg 112 sprays
Applicant: MSD K.K. Approval date: 2012-05-25. Type: Partial change. Active ingredient(s): Mometasone furoate hydrate.
Notes: Drugs with a new additional pediatric dosage for the treatment of allergic rhinitis.